PMDA approval: Avigan Tablet 200 mg
Applicant: Toyama Chemical Co., Ltd. Approval date: 2014-03-24. Type: New approval. Active ingredient(s): Favipiravir.
Notes: A drug with a new active ingredient indicated for novel or re-emerging influenza virus infection s (for use only in patients who have not responded or not responded sufficiently to other anti-influenza virus drugs). [Priority review]